PMDA approval: Votrient Tablets 200 mg
Applicant: GlaxoSmithKline K.K. Approval date: 2012-09-28. Type: New approval. Active ingredient(s): Pazopanib hydrochloride.
Notes: A drug with a new active ingredient indicated for the treatment of soft tissue sarcoma. [Orphan drug]